PMDA approval: Spikevax Intramuscular Injection
Applicant: Moderna Japan Co., Ltd. Approval date: 2023-08-02. Type: Partial change. Active ingredient(s): Elasomeran, Elasomeran and Imelasomeran, Elasomeran and Davesomeran.
Notes: A drug with a new additional pediatric dosage indicated for the prevention of disease caused by SARS-CoV-2 infection (COVID-19). [Drug pertaining to Special Approval for Emergency]